PMDA approval: Finibax for Intravenous Infusion 0.25 g Finibax for Intravenous Infusion 0.5 g Finibax Kit for Intravenous Infusion 0.25 g
Applicant: Shionogi & Co., Ltd. Approval date: 2012-05-25. Type: Partial change. Active ingredient(s): Doripenem hydrate.
Notes: Drugs with a new additional pediatric dosage and with a new additional indication for the treatment of purulent meningitis.